PMDA approval: Vasolan for Intravenous Injection 5 mg
Applicant: Eisai Co., Ltd. Approval date: 2011-05-20. Type: Partial change. Active ingredient(s): Verapamil hydrochloride.
Notes: A drug with a new additional dosage indicated for the treatment of tachyarrhythmia in pediatric patients (paroxysmal supraventricular tachycardia, paroxysmal atrial fibrillation, paroxysmal atrial flutter). [Public knowledge-based application after PAFSC's preliminary assessment]